PMDA approval: Levemir 300 Levemir 300 FlexPen
Applicant: Novo Nordisk Pharma Ltd. Approval date: 2007-10-19. Type: New approval. Active ingredient(s): Insulin detemir (genetical recombination).
Notes: Drugs containing a new active ingredient indicated for the treatment of diabetes mellitus where insulin therapy is indicated.